PMDA approval: (1) Xeloda Tablet 300 (Chugai Pharmaceutical Co., Ltd.) (2) Elplat I.V. Infusion Solution 50 mg (3) Elplat I.V. Infusion Solution 100 mg (4) Elplat I.V. Infusion Solution 200 mg
Applicant: Yakult Honsha Co., Ltd. Approval date: 2015-11-20. Type: Partial change. Active ingredient(s): (1) Capecitabine (2) - (4) Oxaliplatin.
Notes: Drugs with a revised indication for the treatment of gastric cancer.